PMDA approval: Foscavir Infusion Solution 24 mg/mL
Applicant: Clinigen K.K. Approval date: 2019-03-26. Type: Partial change. Active ingredient(s): Foscarnet sodium hydrate.
Notes: A drug with a new additional indication and a new dosage indicated for the treatment of human herpesvirus 6 encephalitis after hematopoietic stem cell transplantation. [Public knowledge-based application after PAFSC's preliminary assessment]